PMDA approval: Rybrevant Intravenous Infusion 350 mg
Applicant: Janssen Pharmaceutical K.K. Approval date: 2025-03-27. Type: Partial change. Active ingredient(s): Amivantamab (genetical recombination).
Notes: A drug with a new indication and a new dosage for the treatment of unresectable advanced or recurrent epidermal growth factor receptor (EGFR) mutation-positive non-small cell lung cancer.